PMDA approval: Allergen Extract for Subcutaneous Injection-HDM "TORII"100,000 JAU/mL Allergen Extract for Subcutaneous Injection-HDM "TORII"10,000 JAU/mL
Applicant: Torii Pharmaceutical Co., Ltd. Approval date: 2014-12-26. Type: New approval. Active ingredient(s): Dermatophagoides farinae extract 10,000 AU/mL, Dermatophagoides pteronyssinus extract 10,000 AU/mL.
Notes: Drugs with new active ingredients indicated for the allergen immunotherapy for house dust mite antigen-induced allergic rhinitis and bronchial asthma.